PMDA approval: Imbruvica Capsules 140 mg
Applicant: Janssen Pharmaceutical K.K. Approval date: 2016-12-02. Type: Partial change. Active ingredient(s): Ibrutinib.
Notes: A drug with a new additional indication and a new dosage for the treatment of relapsed or refractory mantle cell lymphoma. [Orphan drug]